PMDA approval: Campto 40 mg for I.V. infusion Campto 100 mg for I.V. infusion (Yakult Honsha Co., Ltd.) Topotecin Intravenous Drip Infusion 40 mg Topotecin Intravenous Drip Infusion 100 mg (Daiichi Sankyo Company, Limited) Irinotecan Hydrochloride Intravenous Drip Infusion 40 mg "Sawai" Irinotecan Hydrochloride Intravenous Drip Infusion 100 mg "Sawai" (Sawai Pharmaceutical Co., Ltd.) Irinotecan Hydrochloride I.V. Infusion 40 mg "Taiho" Irinotecan Hydrochloride I.V. Infusion 100 mg "Taiho" (Taiho Pharmaceutical Co., Ltd.) Irinotecan Hydrochloride I.V. Infusion 40 mg "Hospira" Irinotecan Hydrochloride I.V. Infusion 100 mg "Hospira"
Applicant: Hospira Japan Co., Ltd. Approval date: 2013-03-25. Type: Partial change. Active ingredient(s): Irinotecan hydrochloride hydrate.
Notes: Drugs with a new additional indication and a new dosage for the treatment of pediatric malignant solid tumor. [Public knowledge-based application after PAFSC's preliminary assessment]